PMDA approval: Nucala for s.c. Injection 100 mg
Applicant: GlaxoSmithKline K.K. Approval date: 2018-05-25. Type: Partial change. Active ingredient(s): Mepolizumab (genetical recombination).
Notes: A drug with a new additional indication and a new dosage for the treatment of eosinophilic granulomatosis with polyangiitis in patients who have not responded sufficiently to conventional treatments. [Orphan drug]